PMDA approval: MaQaid Intravitreal Injection 40 mg
Applicant: Wakamoto Co., Ltd. Approval date: 2017-03-02. Type: Partial change. Active ingredient(s): Triamcinolone acetonide.
Notes: A drug with a new route of administration indicated for alleviation of diabetic macular edema and macular edema associated with retinal vein occlusion or non-infectious uveitis.